PMDA approval: Adcetris for Intravenous Drip Infusion 50 mg
Applicant: Takeda Pharmaceutical Company Limited. Approval date: 2022-05-26. Type: Partial change. Active ingredient(s): Brentuximab vedotin (genetical recombination).
Notes: A drug with a new additional pediatric dosage indicated for the treatment of CD30-positive Hodgkin's lymphoma. [Orphan drug]